PMDA approval: Sumithrin Lotion 5%
Applicant: Kracie Pharma, Ltd. Approval date: 2014-03-24. Type: New approval. Active ingredient(s): Phenothrin.
Notes: A drug with a new indication and a new dosage for the treatment of scabies. [Priority review]